PMDA approval: (1) Tremfya Intravenous Infusion 200 mg (2) Tremfya Subcutaneous Injection 200 mg Syringe (3) Tremfya Subcutaneous Injection 200 mg Pen (4) Tremfya Subcutaneous Injection 100 mg Syringe
Applicant: Janssen Pharmaceutical K.K. Approval date: 2025-03-27. Type: New approval. Active ingredient(s): Guselkumab (genetical recombination).
Notes: (1) A drug with a new route of administration indicated for the remission induction therapy for moderate to severe ulcerative colitis (for use only in patients who have not sufficiently responded to conventional treatments). (2)(3) Drugs with a new indication and a new dosage in an additional dosage form for the maintenance therapy for moderate to severe ulcerative colitis (for use only in patients who have not sufficiently responded to conventional treatments). (4) A drug with a new indication and a new dosage for the maintenance therapy for moderate to severe ulcerative colitis (for use only in patients who have not sufficiently responded to conventional treatments).